PMDA approval: Inavir Dry Powder Inhaler 20 mg
Applicant: Daiichi Sankyo Company, Limited. Approval date: 2013-12-20. Type: Partial change. Active ingredient(s): Laninamivir octanoate hydrate.
Notes: A drug with a new additional indication and a new dosage for prophylaxis of influenza A or B virus infections.